PMDA approval: Venclexta Tablets 10 mg Venclexta Tablets 50 mg Venclexta Tablets 100 mg
Applicant: AbbVie GK. Approval date: 2025-03-27. Type: Partial change. Active ingredient(s): Venetoclax.
Notes: Drugs with a new indication and a new dosage for the treatment of relapsed or refractory mantle cell lymphoma. [Orphan drug]